PMDA approval: Kidparen Injection
Applicant: Otsuka Pharmaceutical Factory, Inc. Approval date: 2024-09-24. Type: New approval. Active ingredient(s): N/A for this combination drug.
Notes: A combination prescription drug with similar formulations indicated for the supplementation of water, electrolytes, calories, amino acids, and vitamins in patients with chronic renal failure (for use only in patients with or at a risk of hyperkalemia or hyperphosphatemia) for whom receiving oral or enteral nutrition is impossible or insufficient and the total parenteral nutrition is the only choice.